PMDA approval: Vizamyl Intravenous Injectable
Applicant: Nihon Medi-Physics Co., Ltd. Approval date: 2017-09-27. Type: New approval. Active ingredient(s): Flutemetamol (18F).
Notes: A drug with a new active ingredient indicated for visualization of beta ‑amyloid plaques in the brains of patients with cognitive impairment suspected to be Alzheimer's disease.